PMDA approval: L'estrogel 0.06%
Applicant: Shiseido Company, Limited. Approval date: 2011-11-25. Type: Partial change. Active ingredient(s): Estradiol.
Notes: A drug with a new dosage. This drug is indicated for the treatment of vasomotor symptoms (hot flush and sweating) associated with menopausal disorder and ovarian deficiency symptoms.